PMDA approval: Tevimbra I.V. Infusion 100 mg
Applicant: BeiGene Japan GK. Approval date: 2025-03-27. Type: New approval. Active ingredient(s): Tislelizumab (genetical recombination).
Notes: A drug with a new active ingredient indicated for the treatment of unresectable advanced or recurrent esophageal cancer.